PMDA approval: Humira 40 mg for S.C. Injection Syringe 0.8 mL
Applicant: Abbvie G.K. Approval date: 2013-05-16. Type: Partial change. Active ingredient(s): Adalimumab (genetical recombination).
Notes: A drug with a new additional indication and a new dosage for the treatment of intestinal Behcet's disease in patients who have not responded sufficiently to conventional treatments.